PMDA approval: Pomalyst Capsules 1 mg Pomalyst Capsules 2 mg Pomalyst Capsules 3 mg Pomalyst Capsules 4 mg
Applicant: Celgene K.K. Approval date: 2015-03-26. Type: New approval. Active ingredient(s): Pomalidomide.
Notes: Drugs with a new active ingredient indicated for the treatment of relapsed or refractory multiple myeloma. [Orphan drug]